PMDA approval: Parmodia Tab. 0.1 mg
Applicant: Kowa Company, Ltd. Approval date: 2017-07-03. Type: New approval. Active ingredient(s): Pemafibrate.
Notes: A drug with a new active ingredient indicated for the treatment of hyperlipidaemia (including familial hyperlipidaemia).